PMDA approval: Revlimid Capsules 5 mg
Applicant: Celgene K.K. Approval date: 2010-06-25. Type: New approval. Active ingredient(s): Lenalidomide hydrate.
Notes: A drug with a new active ingredient indicated for the treatment of relapsed or refractory multiple myeloma. [Orphan drug]